PMDA approval: Lympack TA5
Applicant: Nipro Corporation. Approval date: 2022-03-28. Type: New approval. Active ingredient(s): N/A for this combination drug.
Notes: A combination prescription drug with similar formulations to be used as perfusion fluids for hemodialysis in patients with chronic renal failure (use in any of the following cases: ○ when bicarbonate dialysate with high bicarbonate concentration may cause excessive alkalosis; ○ when the patient’s blood sugar level is difficult to control with sugarless dialysate; ○ when other bicarbonate dialysate is not sufficiently effective to improve hyperkalemia and hypermagnesemia or may cause hypercalcemia).